PMDA approval: Micamlo Combination Tablets AP
Applicant: Nippon Boehringer Ingelheim Co., Ltd. Approval date: 2010-07-23. Type: New approval. Active ingredient(s): Telmisartan/amlodipine besilate.
Notes: A new combination drug indicated for the treatment of hypertension.